PMDA approval: Copegus Tablet 200 mg
Applicant: Chugai Pharmaceutical Co., Ltd. Approval date: 2017-03-24. Type: Partial change. Active ingredient(s): Ribavirin.
Notes: A drug with a new additional indication for the improvement of viraemia in patients with chronic hepatitis C or compensated cirrhosis type C in neither Serogroup 1 (genotype 1) nor Serogroup 2 (genotype 2). [Expedited review]